PMDA approval: (1) Isovorin Injection 25 mg Isovorin Injection 100 mg (Pfizer Japan Inc.) (2) Levofolinate for I.V. Drip Infusion 25 "Ohara" Levofolinate for I.V. Drip Infusion 100 "Ohara" (Ohara Pharmaceutical Co., Ltd.) (3) Levofolinate for I.V. Infusion 25 mg “Yakult” Levofolinate for I.V. Infusion 100 mg “Yakult” (Yakult Honsha Co., Ltd.) (4) Levofolinate for I.V. Infusion 25 mg "NK" Levofolinate for I.V. Infusion100 mg "NK" (Takata Pharmaceutical Co.,Ltd.) (5) Levofolinate for I.V. Infusion 25 mg "NP" Levofolinate for I.V. Infusion 100 mg "NP"
Applicant: Nipro Corporation. Approval date: 2018-09-21. Type: Partial change. Active ingredient(s): Except (2): Levofolinate calcium (2): Calcium levofolinate hydrate.
Notes: Drugs with a new indication and a new dosage for the treatment of small intestine cancer. [Public knowledge-based application after PAFSC's preliminary assessment]